PMDA approval: Tecvayli Subcutaneous Injection 153 mg Tecvayli Subcutaneous Injection 30 mg
Applicant: Janssen Pharmaceutical K.K. Approval date: 2024-12-27. Type: New approval. Active ingredient(s): Teclistamab (genetical recombination).
Notes: Drugs with a new active ingredient indicated for the treatment of relapsed or refractory multiple myeloma (for use only if refractory or intolerant to standard therapies). [Orphan drug]